PMDA approval: Amlodipine Tab. 2.5 mg "Amel" Amlodipine Tab. 5 mg "Amel" Amlodipine OD Tab. 2.5 mg "Amel" Amlodipine OD Tab. 5 mg "Amel"
Applicant: Kyowa Pharmaceutical Industry Co., Ltd. Approval date: 2012-06-22. Type: Partial change. Active ingredient(s): Amlodipine besilate.
Notes: Drugs with a new additional pediatric dosages. These drugs are indicated for the treatment of hypertension. [Public knowledge-based application after PAFSC's preliminary assessment]